PMDA approval: Sogroya Subcutaneous Injection 5 mg Sogroya Subcutaneous Injection 10 mg
Applicant: Novo Nordisk Pharma Ltd. Approval date: 2021-01-22. Type: New approval. Active ingredient(s): Somapacitan (genetical recombination).
Notes: Drugs with a new active ingredient indicated for the treatment of adult growth hormone deficiency (used only for severe cases).